PMDA approval: Galafold Capsules 123 mg
Applicant: Amicus Therapeutics, Inc. Approval date: 2022-12-23. Type: Partial change. Active ingredient(s): Migalastat hydrochloride.
Notes: A drug with a new additional pediatric dosage indicated for the treatment of Fabry disease in patients with GLA mutations categorized as amenable to treatment with migalastat. [Orphan drug]